PMDA approval: Epadel Capsules 300 Epadel S300 Epadel S600 Epadel S900
Applicant: Mochida Pharmaceutical Co., Ltd. Approval date: 2012-06-22. Type: Partial change. Active ingredient(s): Ethyl icosapentate.
Notes: Drugs with a new dosage. These drugs are indicated for the treatment of hyperlipemia.